PMDA approval: Actonel Tablets 17.5 mg (Takeda Pharmacdeutical Co., Ltd.) Benet Tablets 17.5 mg
Applicant: Ajinomoto Co., Inc. Approval date: 2007-04-18. Type: Partial change. Active ingredient(s): Risedronate sodium hydrate.
Notes: Drugs with a new dosage and in a new dosage form indicated for the treatment of osteoporosis (once-weekly dosing).